PMDA approval: Ancaron Inj. 150
Applicant: Sanofi K.K. Approval date: 2013-05-31. Type: Partial change. Active ingredient(s): Amiodarone hydrochloride.
Notes: A drug with a new additional indication and a new dosage indicated for the treatment of cardiac arrest due to ventricular fibrillation/pulseless ventricular tachycardia, resistant to electrical cardioversion.